PMDA approval: Zacras Combination Tablets LD Zacras Combination Tablets HD
Applicant: Takeda Pharmaceutical Company Limited. Approval date: 2014-03-24. Type: New approval. Active ingredient(s): Azilsartan/amlodipine besilate.
Notes: New combination drugs indicated for the treatment of hypertension.